PMDA approval: Zavicefta Combination for Intravenous Infusion
Applicant: Pfizer Japan Inc. Approval date: 2024-06-24. Type: New approval. Active ingredient(s): Avibactam sodium/Ceftazidime hydrate.
Notes: A new combination drug with a new active ingredient indicated for the treatment of sepsis, pneumonia, cystitis, pyelonephritis, peritonitis, intra-abdominal abscess, cholecystitis, and liver abscess caused by Zavicefta-sensitive Escherichia coli, Citrobacter spp., Klebsiella spp., Enterobacter spp., Serratia spp., Proteus spp., Morganella morganii, Haemophilus influenzae, and Pseudomonas aeruginosa.